PMDA approval: Padcev for I.V. infusion 30 mg
Applicant: Astellas Pharma Inc. Approval date: 2021-09-27. Type: New approval. Active ingredient(s): Enfortumab vedotin (genetical recombination).
Notes: A drug with a new active ingredient indicated for the treatment of unresectable urothelial carcinoma that has progressed after cancer chemotherapy. [Priority review]